PMDA approval: Epclusa Combination Tablets
Applicant: Gilead Sciences K.K. Approval date: 2019-01-08. Type: New approval. Active ingredient(s): Sofosbuvir/Velpatasvir.
Notes: A new combination drug with a new active ingredient indicated for: - Improvement of viremia in patients with chronic hepatitis C or compensated cirrhosis type C who have previously been treated. - Improvement of viremia in patients with decompensated cirrhosis type C. [Priority review]